PMDA approval: Zilbrysq Syringe for S.C. Injections 16.6 mg Zilbrysq Syringe for S.C. Injections 23.0 mg Zilbrysq Syringe for S.C. Injections 32.4 mg
Applicant: UCB Japan Co., Ltd. Approval date: 2023-09-25. Type: New approval. Active ingredient(s): Zilucoplan sodium.
Notes: Drugs with a new active ingredient indicated for the treatment of generalized myasthenia gravis (for use only in patients who have not sufficiently responded to steroids or other immunosuppressants).